PMDA approval: Alecensa Capsule 150 mg
Applicant: Chugai Pharmaceutical Co., Ltd. Approval date: 2020-02-21. Type: Partial change. Active ingredient(s): Alectinib hydrochloride.
Notes: A drug with a new indication and a new dosage for the treatment of relapsed or refractory ALK fusion gene-positive anaplastic large cell lymphoma. [Orphan drug]